PMDA approval: Metastron Injectable
Applicant: GE Healthcare Ltd. Approval date: 2007-07-31. Type: New approval. Active ingredient(s): Strontium (89Sr) chloride.
Notes: A drug containing a new active ingredient indicated for pain relief in bone scintigraphy-positive metastatic lesions in patients with solid cancers.